PMDA approval: Pegasys S.C. 90 μg Copegus Tablets 200 mg
Applicant: Chugai Pharmaceutical Co., Ltd. Approval date: 2011-07-01. Type: Partial change. Active ingredient(s): Peginterferon alfa-2a (genetical recombination) Ribavirin.
Notes: Drugs with a new additional indication for the improvement of viremia in patients with compensated cirrhosis C. A new dosage of Pegasis S.C. 90 μg has also been approved. [Priority review]